PMDA approval: Tasigna Capsules 50 mg Tasigna Capsules 150 mg Tasigna Capsules 200 mg
Applicant: Novartis Pharma K.K. Approval date: 2017-12-25. Type: Partial change. Active ingredient(s): Nilotinib hydrochloride hydrate.
Notes: Drugs with a new pediatric dosage indicated for the treatment of chronic- or accelerated-phase chronic myeloid leukemia. [Expedited review (only Tasigna Capsules 50 mg)]